PMDA approval: Trazenta Tablets 5 mg
Applicant: Nippon Boehringer Ingelheim Co., Ltd. Approval date: 2013-03-25. Type: Partial change. Active ingredient(s): Linagliptin.
Notes: A drug with a new indication for the treatment of type 2 diabetes mellitus.